PMDA approval: Keytruda Injection 100 mg
Applicant: MSD K.K. Approval date: 2025-05-19. Type: Partial change. Active ingredient(s): Pembrolizumab (genetical recombination).
Notes: A drug with a new indication for the treatment of unresectable advanced or recurrent malignant pleural mesothelioma.